PMDA approval: Actonel Tab. 17.5 mg (Ajinomoto Co., Inc.) Benet Tablets 17.5 mg.
Applicant: Takeda Pharmacdeutical Co., Ltd. Approval date: 2008-07-16. Type: Partial change. Active ingredient(s): Risedronate sodium hydrate.
Notes: Drugs with a new indication and a new dosage for the treatment of Paget's disease of bone. [Orphan drug]